PMDA approval: Mayzent Tablets 0.25 mg Mayzent Tablets 2 mg
Applicant: Novartis Pharma K.K. Approval date: 2020-06-29. Type: New approval. Active ingredient(s): Siponimod fumaric acid.
Notes: Drugs with a new active ingredient indicated for the prevention of relapse and for delaying the accumulation of physical disability in secondary progressive multiple sclerosis (SPMS). [Orphan drug]